PMDA approval: Vyxeos Combination for I.V. Injection
Applicant: Nippon Shinyaku Co., Ltd. Approval date: 2024-03-26. Type: New approval. Active ingredient(s): Daunorubicin hydrochloride, Cytarabine.
Notes: A new combination drug indicated for the treatment of high-risk acute myeloid leukemia. [Orphan drug]